PMDA approval: Kavigale Injection Solution 300 mg
Applicant: AstraZeneca K.K. Approval date: 2024-12-27. Type: New approval. Active ingredient(s): Sipavibart (genetical recombination).
Notes: A drug with a new active ingredient indicated for the prevention of disease caused by SARS-CoV-2 infection (COVID-19). [Expedited review]